PMDA approval: Aldreb for Injection 150 mg
Applicant: GlaxoSmithKline K.K. Approval date: 2015-03-26. Type: New approval. Active ingredient(s): Colistin sodium methanesulfonate.
Notes: A drug with a new route of administration indicated for the treatment of infections caused by colistin-sensitive Escherichia coli, Citrobacter, Klebsiella, Enterobacter, Pseudomonas aeruginosa, and Acinetobacter (limited to the strains resistant to other antimicrobial drugs). [Orphan drug]